PMDA approval: Brineura Intracerebroventricular Injectable Solution 150 mg
Applicant: BioMarin Pharmaceutical Japan K.K. Approval date: 2019-09-20. Type: New approval. Active ingredient(s): Cerliponase alfa (genetical recombination).
Notes: A drug with a new active ingredient indicated for the treatment of neuronal ceroid lipofuscinosis type 2 (CLN2). [Orphan drug]